PMDA approval: Spikevax Intramuscular Injection
Applicant: Moderna Japan Co., Ltd. Approval date: 2023-10-24. Type: Partial change. Active ingredient(s): Elasomeran, Elasomeran and Imelasomeran, Elasomeran and Davesomeran, Andusomeran.
Notes: A drug with a new dosage for the prevention of disease caused by SARS-CoV-2 infection (COVID- 19). [Drug pertaining to Special Approval for Emergency]